PMDA approval: Ibulief I.V. Injection 20 mg
Applicant: Senju Pharmaceutical Co., Ltd. Approval date: 2018-01-19. Type: New approval. Active ingredient(s): Ibuprofen L-lysine.
Notes: A drug with a new active ingredient indicated for the treatment of patent ductus arteriosus in premature infants when conservative treatment (fluid restriction, diuretic use, etc.) is not sufficiently effective.